PMDA approval: Nexavar Tablets 200 mg
Applicant: Bayer Yakuhin, Ltd. Approval date: 2016-02-29. Type: Partial change. Active ingredient(s): Sorafenib tosilate.
Notes: A drug with a revised indication for the treatment of unresectable thyroid cancer. [Orphan drug]